PMDA approval: Ozempic Subcutaneous Injection 2 mg
Applicant: Novo Nordisk Pharma Ltd. Approval date: 2018-03-23. Type: New approval. Active ingredient(s): Semaglutide (genetical recombination).
Notes: A drug with a new active ingredient indicated for the treatment of type 2 diabetes mellitus.